PMDA approval: Hemlibra s.c. 30 mg Hemlibra s.c. 60 mg Hemlibra s.c. 90 mg Hemlibra s.c. 105 mg Hemlibra s.c. 150 mg
Applicant: Chugai Pharmaceutical Co., Ltd. Approval date: 2022-06-20. Type: Partial change. Active ingredient(s): Emicizumab (genetical recombination).
Notes: Drugs with a new indication and a new dosage for the control of bleeding tendency in patients with acquired hemophilia A. [Orphan drug]